PMDA approval: Buccolam Oromucosal Solution 2.5 mg Buccolam Oromucosal Solution 5 mg Buccolam Oromucosal Solution 7.5 mg Buccolam Oromucosal Solution 10 mg
Applicant: Takeda Pharmaceutical Company Limited. Approval date: 2020-09-25. Type: New approval. Active ingredient(s): Midazolam.
Notes: Drugs with a new route of administration indicated for the treatment of status epilepticus. [Orphan drug]